PMDA approval: Efient Tablets 3.75 mg Efient Tablets 5 mg
Applicant: Daiichi Sankyo Company, Limited. Approval date: 2014-03-24. Type: New approval. Active ingredient(s): Prasugrel hydrochloride.
Notes: Drugs with a new active ingredient indicated for the treatment of the following ischemic heart diseases managed with percutaneous coronary intervention (PCI): acute coronary syndrome (unstable angina, non ST-segment elevation myocardial infarction, ST-segment elevation myocardial infarction), stable angina, old myocardial infarction.